PMDA approval: Pifeltro Tablets 100 mg
Applicant: MSD K.K. Approval date: 2020-01-14. Type: New approval. Active ingredient(s): Doravirine.
Notes: A drug with a new active ingredient indicated for the treatment of HIV-1 infection. [Orphan drug]